PMDA approval: EOB・Primovist Injection Syringe
Applicant: Bayer Yakuhin Ltd. Approval date: 2007-10-19. Type: New approval. Active ingredient(s): Gadoxetate sodium.
Notes: A drug containing a new active ingredient indicated for use in magnetic resonance computerized tomography imaging of liver tumors.